PMDA approval: Eylea Intravitreal Injection 40 mg/mL Eylea Intravitreal Injection Kit 40 mg/mL
Applicant: Bayer Yakuhin, Ltd. Approval date: 2014-11-18. Type: Partial change. Active ingredient(s): Aflibercept (genetical recombination).
Notes: Drugs with a new additional indication and a new dosage for the treatment of diabetic macular edema.